PMDA approval: Zestril Tablets 5 Zestril Tablets 10 Zestril Tablets 20
Applicant: AstraZeneca K.K. Approval date: 2012-06-22. Type: Partial change. Active ingredient(s): Lisinopril hydrate.
Notes: Drugs with a new additional pediatric dosage. These drugs are indicated for the treatment of hypertension. [Public knowledge-based application after PAFSC's preliminary assessment]